PMDA approval: Tafinlar Capsules 50 mg Tafinlar Capsules 75 mg
Applicant: Novartis Pharma K.K. Approval date: 2016-03-28. Type: New approval. Active ingredient(s): Dabrafenib mesilate.
Notes: Drugs with a new active ingredient indicated for the treatment of unresectable melanoma with BRAF mutation. [Orphan drug]